PMDA approval: Nucala for s.c. Injection 100 mg
Applicant: GlaxoSmithKline K.K. Approval date: 2016-03-28. Type: New approval. Active ingredient(s): Mepolizumab (genetical recombination).
Notes: A drug with a new active ingredient indicated for the treatment of bronchial asthma (for use only in patients with intractable bronchial asthma whose asthmatic responses are uncontrollable with conventional therapies).